PMDA approval: Korsuva IV Injection Syringe for Dialysis 17.5 µg Korsuva IV Injection Syringe for Dialysis 25.0 µg Korsuva IV Injection Syringe for Dialysis 35.0 µg
Applicant: Maruishi Pharmaceutical Co., Ltd. Approval date: 2023-09-25. Type: New approval. Active ingredient(s): Difelikefalin acetate.
Notes: Drugs with a new active ingredient indicated for the improvement of pruritus in patients on hemodialysis (for use only in patients who have not sufficiently responded to conventional treatments).